PMDA approval: Sulprep Combination Solution
Applicant: Nihon Pharmaceutical Co., Ltd. Approval date: 2021-01-22. Type: New approval. Active ingredient(s): Anhydrous sodium sulfate, Potassium sulfate, Magnesium sulfate hydrate.
Notes: A new combination drug indicated for bowel cleansing preparation for colonoscopy.